PMDA approval: DaTscan Intravenous Injection
Applicant: Nihon Medi-Physics Co., Ltd. Approval date: 2013-09-20. Type: New approval. Active ingredient(s): Ioflupane (123I).
Notes: A drug with a new active ingredient indicated for dopamine transporter scintigraphy in the diagnoses of Parkinson's syndrome and dementia with Lewy bodies.